PMDA approval: Lixiana Tablets 15 mg Lixiana Tablets 30 mg
Applicant: Daiichi Sankyo Company, Limited. Approval date: 2011-04-22. Type: New approval. Active ingredient(s): Edoxaban tosilate hydrate.
Notes: Drugs with a new active ingredient indicated for prevention of venous thromboembolism in patients undergoing orthopedic surgery of lower limbs including total knee arthroplasty, total hip arthroplasty and hip fracture surgery.